PMDA approval: Iressa Tablets 250
Applicant: AstraZeneca K.K. Approval date: 2011-11-25. Type: Partial change. Active ingredient(s): Gefitinib.
Notes: A drug with a revised indication for the treatment of unresectable advanced or recurrent non-small cell lung cancer in patients with EGFR gene mutation.